PMDA approval: Signifor LAR Kit for i.m. injection 20 mg Signifor LAR Kit for i.m. injection 40 mg Signifor LAR Kit for i.m. injection 60 mg
Applicant: Novartis Pharma K.K. Approval date: 2016-09-28. Type: New approval. Active ingredient(s): Pasireotide pamoate.
Notes: Drugs with a new active ingredient indicated for the improvement of hypersecretion of growth hormone and IGF-I (somatomedin-C) and related symptoms in acromegaly and pituitary gigantism (when surgical therapies are not sufficiently effective or are difficult to perform).